PMDA approval: Afstyla I.V. Injection 250 Afstyla I.V. Injection 500 Afstyla I.V. Injection 1000 Afstyla I.V. Injection 1500 Afstyla I.V. Injection 2000 Afstyla I.V. Injection 2500 Afstyla I.V. Injection 3000
Applicant: CSL Behring K.K. Approval date: 2017-09-27. Type: New approval. Active ingredient(s): Lonoctocog alfa (genetical recombination).
Notes: Drugs with a new active ingredient indicated for the control of bleeding tendency in patients with blood coagulation factor VIII deficiency.